PMDA approval: Samtirel Oral Suspension 15%
Applicant: GlaxoSmithKline K.K. Approval date: 2012-01-18. Type: New approval. Active ingredient(s): Atovaquone.
Notes: A drug with a new active ingredient indicated for the treatment and prevention of Pneumocystis pneumonia. [Priority review]